PMDA approval: ColomforT Oral Suspension 25%
Applicant: Fushimi Pharmaceutical Co.,Ltd. Approval date: 2016-03-28. Type: New approval. Active ingredient(s): Barium sulfate.
Notes: A drug with a new indication and a new dosage in an additional dosage form indicated for the support of computed tomographic colonography with labelling of bowel contents.